PMDA approval: Loqoa Tape
Applicant: Taisho Pharmaceutical Co., Ltd. Approval date: 2015-09-28. Type: New approval. Active ingredient(s): Esflurbiprofen/Mentha oil.
Notes: A new combination drug with a new active ingredient indicated for the relief of inflammation and pain associated with osteoarthritis.